PMDA approval: Romiplate for S.C. Injection 250 µg
Applicant: Kyowa Kirin Co., Ltd. Approval date: 2023-09-25. Type: Partial change. Active ingredient(s): Romiplostim (genetical recombination).
Notes: A drug with a new indication and a new dosage for the treatment of aplastic anemia.